PMDA approval: Unituxin I.V. Injection 17.5 mg/5 mL
Applicant: Ohara Pharmaceutical Co.,Ltd. Approval date: 2021-06-23. Type: New approval. Active ingredient(s): Dinutuximab (genetical recombination).
Notes: A drug with a new active ingredient indicated for the treatment of neuroblastoma following high-dose chemotherapy. [Orphan drug]